PMDA approval: Orkedia Tablets 1 mg Orkedia Tablets 2 mg
Applicant: Kyowa Hakko Kirin Co., Ltd. Approval date: 2018-03-23. Type: New approval. Active ingredient(s): Evocalcet.
Notes: Drugs with a new active ingredient indicated for the treatment of secondary hyperparathyroidism in patients on maintenance dialysis.